PMDA approval: OxyFast Injection 10 mg OxyFast Injection 50 mg
Applicant: Shionogi & Co., Ltd. Approval date: 2012-01-18. Type: New approval. Active ingredient(s): Oxycodone hydrochloride hydrate.
Notes: Drugs with a new route of administration indicated for analgesia in various types of cancer with moderate to severe pain.